PMDA approval: Lenvima Capsule 4 mg
Applicant: Eisai Co., Ltd. Approval date: 2018-03-23. Type: Partial change. Active ingredient(s): Lenvatinib mesilate.
Notes: A drug with a new additional indication and a new dosage indicated for the treatment of unresectable hepatic cell carcinoma.